PMDA approval: Orapenem Fine Granules 10% for Pediatric
Applicant: Meiji Seika Kaisha, Ltd. Approval date: 2009-04-22. Type: New approval. Active ingredient(s): Tebipenem pivoxil.
Notes: A drug with a new active ingredient indicated for the treatment of pneumonia, otitis media, and sinusitis.